PMDA approval: Truvada Combination Tab.
Applicant: Gilead Sciences, Inc. Approval date: 2024-08-28. Type: Partial change. Active ingredient(s): Emtricitabine/ Tenofovir disoproxil fumarate.
Notes: A drug with a new indication and a new dosage for the pre-exposure prophylaxis against HIV-1 infection. [Public knowledge-based application after PAFSC's preliminary assessment]